PMDA approval: Lixiana Tablets 15 mg Lixiana OD Tablets 15 mg
Applicant: Daiichi Sankyo Company, Limited. Approval date: 2021-08-25. Type: Partial change. Active ingredient(s): Edoxaban tosilate hydrate.
Notes: Drugs with a new dosage indicated for the prevention of ischemic stroke and systemic embolism in patients with non-valvular atrial fibrillation.